PMDA approval: Dupixent 300 mg Syringe for S.C. Injection
Applicant: Sanofi K.K. Approval date: 2019-03-26. Type: Partial change. Active ingredient(s): Dupilumab (genetical recombination).
Notes: A drug with a new additional indication, a new dosage and other characteristics for the treatment of bronchial asthma (for use only in patients with severe or intractable bronchial asthma whose asthmatic responses are uncontrollable with conventional therapies).